PMDA approval: Tecfidera Capsules 120 mg Tecfidera Capsules 240 mg
Applicant: Biogen Japan Ltd. Approval date: 2016-12-19. Type: New approval. Active ingredient(s): Dimethyl fumarate.
Notes: Drugs with a new active ingredient indicated for the prevention of relapse and for delaying the accumulation of physical disability in multiple sclerosis. [Orphan drug]